PMDA approval: Xocova Tablets 125 mg
Applicant: Shionogi & Co., Ltd. Approval date: 2022-11-22. Type: New approval. Active ingredient(s): Ensitrelvir fumaric acid.
Notes: A drug with a new active ingredient indicated for the treatment of disease caused by SARS-CoV-2 infection (COVID-19). [Drug pertaining to Emergency Approval]